PMDA approval: Eylea Intravitreal Injection 40 mg/mL, Eylea Intravitreal Injection Kit 40 mg/mL
Applicant: Bayer Yakuhin, Ltd. Approval date: 2012-09-28. Type: New approval. Active ingredient(s): Aflibercept (genetical recombination).
Notes: Drugs with a new active ingredient indicated for the treatment of age-related macular degeneration associated with subfoveal choroidal neovascularization.